PMDA approval: Lixiana Tablets 15 mg Lixiana Tablets 30 mg Lixiana Tablets 60 mg
Applicant: Daiichi Sankyo Company, Limited. Approval date: 2014-09-26. Type: Partial change. Active ingredient(s): Edoxaban tosilate hydrate.
Notes: Drugs with new additional indications and new dosages, and a drug with a newly-added dosage form indicated for prevention of ischemic stroke and systemic embolism in patients with non-valvular atrial fibrillation, or for the treatment and prevention of the recurrence of venous thromboembolism (deep vein thrombosis and pulmonary thromboembolism).